PMDA approval: Pluvicto Injection
Applicant: Novartis Pharma K.K. Approval date: 2025-09-19. Type: New approval. Active ingredient(s): Lutetium (177Lu) vipivotide tetraxetan.
Notes: A drug with a new active ingredient indicated for the treatment of PSMA-positive, metastatic castration-resistant prostate cancer.